PMDA approval: Thyrogen IM Injection 0.9 mg
Applicant: Sato Pharmaceutical Co., Ltd. Approval date: 2008-10-16. Type: New approval. Active ingredient(s): Thyrotropin human alfa (genetical recombination).
Notes: A drug containing a new active ingredient indicated for use as an adjunctive diagnostic tool for serum thyroglobulin (Tg) testing with or without radioiodine scintigraphy in patients with differentiated thyroid cancer who have undergone near total or total thyroidectomy. [Orphan drug]